PMDA approval: Parmodia XR Tablets 0.2 mg Parmodia XR Tablets 0.4 mg
Applicant: Kowa Company, Ltd. Approval date: 2023-06-26. Type: New approval. Active ingredient(s): Pemafibrate.
Notes: Drugs in a new dosage form indicated for the treatment of hyperlipidaemia (including familial hyperlipidaemia).